PMDA approval: Dinagest Tablets 1 mg
Applicant: Mochida Pharmaceutical Co., Ltd. Approval date: 2007-10-19. Type: New approval. Active ingredient(s): Dienogest.
Notes: A drug containing a new active ingredient indicated for the treatment of endometriosis.